PMDA approval: Mucosta Ophthalmic Solution UD 2%
Applicant: Otsuka Pharmaceutical Co., Ltd. Approval date: 2011-09-26. Type: New approval. Active ingredient(s): Rebamipide.
Notes: A drug with a new route of administration indicated for the treatment of dry eye.